PMDA approval: Abrysvo Intramuscular Injection
Applicant: Pfizer Japan Inc. Approval date: 2024-01-18. Type: New approval. Active ingredient(s): Respiratory syncytial virus-A prefusion F protein Respiratory syncytial virus-B prefusion F protein.
Notes: A drug with a new active ingredient indicated for active immunization of pregnant individuals for the prevention of lower respiratory tract disease caused by respiratory syncytial virus in neonates and infants.